PMDA approval: Inavir for Inhalation Suspension 160 mg Set
Applicant: Daiichi Sankyo Company, Limited. Approval date: 2019-06-18. Type: New approval. Active ingredient(s): Laninamivir octanoate hydrate.
Notes: A drug in a new dosage form indicated for the treatment of influenza A or B virus infection.